PMDA approval: Pegintron Powder for Injection 50 μg/0.5 mL Pegintron Powder for Injection 100 μg/0.5 mL Pegintron Powder for Injection 150 μg/0.5 mL
Applicant: MSD K.K. Approval date: 2015-05-26. Type: Partial change. Active ingredient(s): Peginterferon alfa-2b (genetical recombination).
Notes: Drugs with a new additional indication and a new dosage for the adjuvant treatment of melanoma. [Orphan drug]